PMDA approval: Basen Tablets 0.2 Basen OD Tablets 0.2
Applicant: Takeda Pharmaceutical Company Limited. Approval date: 2009-10-16. Type: Partial change. Active ingredient(s): Voglibose.
Notes: Drugs with a new additional indication and a new dosage for prevention of type 2 diabetes mellitus in patients with impaired glucose tolerance (only for whom glycemic control is not sufficient by diet and/or exercise).